PMDA approval: Fycompa for Intravenous Infusion 2 mg
Applicant: Eisai Co., Ltd. Approval date: 2024-01-18. Type: New approval. Active ingredient(s): Perampanel hydrate.
Notes: A drug with a new route of administration indicated for the following treatment: partial seizures (including secondary generalized seizures) in patients with epilepsy and an adjunctive therapy with other antiepileptic drugs to treat tonic-clonic seizure in patients with epilepsy who have not responded sufficiently to other antiepileptic drugs. It is used as an alternative therapy for perampanel oral formulation in patients who are temporarily unable to be administered orally.